PMDA approval: Otezla Tablets 10 mg Otezla Tablets 20 mg Otezla Tablets 30 mg
Applicant: Amgen K.K. Approval date: 2025-03-27. Type: Partial change. Active ingredient(s): Apremilast.
Notes: Drugs with a new indication for the treatment of palmoplantar pustulosis in patients who have not responded sufficiently to local treatment.